PMDA approval: Esperoct for I.V. Injection 500 Esperoct for I.V. Injection 1000 Esperoct for I.V. Injection 1500 Esperoct for I.V. Injection 2000 Esperoct for I.V. Injection 3000
Applicant: Novo Nordisk Pharma Ltd. Approval date: 2019-09-20. Type: New approval. Active ingredient(s): Turoctocog alfa pegol (genetical recombination).
Notes: Drugs with a new active ingredient indicated for the control of bleeding tendency in patients with blood coagulation factor VIII deficiency.